PMDA approval: Zeposia Capsules 0.92 mg Zeposia Capsules Starter Pack
Applicant: Bristol-Myers Squibb K.K. Approval date: 2024-12-27. Type: New approval. Active ingredient(s): Ozanimod hydrochloride.
Notes: Drugs with a new active ingredient indicated for the treatment of moderate to severe ulcerative colitis (for use only in patients who have not sufficiently responded to conventional treatments).